PMDA approval: Minirinmelt OD Tablets 25 μg Minirinmelt OD Tablets 50 μg
Applicant: Ferring Pharmaceuticals Co., Ltd. Approval date: 2019-06-18. Type: New approval. Active ingredient(s): Desmopressin acetate hydrate.
Notes: Drugs with a new additional indication and a new dosage in an additional dosage form indicated for the treatment of nocturia due to nocturnal polyuria in males.